PMDA approval: Veklury for Intravenous Injection 100 mg (Solution) Veklury for Intravenous Injection 100 mg (Lyophilized powder)
Applicant: Gilead Sciences K.K. Approval date: 2020-05-07. Type: New approval. Active ingredient(s): Remdesivir.
Notes: Drugs with a new active ingredient indicated for the treatment of disease caused by SARS-CoV-2 infection (COVID-19). [Drug pertaining to Special Approval for Emergency]